PMDA approval: Revlimid Capsules 2.5 mg Revlimid Capsules 5 mg
Applicant: Celgene K.K. Approval date: 2020-02-21. Type: Partial change. Active ingredient(s): Lenalidomide hydrate.
Notes: Drugs with new indications and a new dosage for the treatment of relapsed or refractory follicular lymphoma and marginal zone lymphoma.